PMDA approval: Rinvoq Tablets 7.5 mg Rinvoq Tablets 15 mg
Applicant: AbbVie GK. Approval date: 2020-01-23. Type: New approval. Active ingredient(s): Upadacitinib hydrate.
Notes: Drugs with a new active ingredient indicated for the treatment of rheumatoid arthritis (including the prevention of structural joint damage) in patients who have not responded sufficiently to conventional treatments.